PMDA approval: Onivyde I.V. Infusion 43 mg
Applicant: Nihon Servier Co., Ltd. Approval date: 2020-03-25. Type: New approval. Active ingredient(s): Irinotecan hydrochloride hydrate.
Notes: A drug with a new indication in a new dosage form for the treatment of unresectable pancreatic cancer that has progressed after cancer chemotherapy.